PMDA approval: Vancomycin Ophthalmic Ointment 1%
Applicant: Toa Pharmaceutical Co., Ltd. Approval date: 2009-10-16. Type: New approval. Active ingredient(s): Vancomycin hydrochloride.
Notes: A drug with a new route of administration indicated for the treatment of conjunctivitis, blepharitis, meibomianitis, and dacryocystitis caused by vancomycin-sensitive methicillin-resistant Staphylococcus aureus (MRSA) and methicillin-resistant Staphylococcus epidermidis (MRSE). [Orphan drug]